PMDA approval: Perjeta Intravenous Infusion 420 mg/14 mL
Applicant: Chugai Pharmaceutical Co., Ltd. Approval date: 2018-10-10. Type: Partial change. Active ingredient(s): Pertuzumab (genetical recombination).
Notes: A drug with a new indication for the treatment of HER2-positive breast cancer.